PMDA approval: Gazyva Intravenous Infusion 1000 mg
Applicant: Chugai Pharmaceutical Co., Ltd. Approval date: 2022-12-23. Type: Partial change. Active ingredient(s): Obinutuzumab (genetical recombination).
Notes: A drug with a new indication and a new dosage for the treatment of CD20-positive chronic lymphocytic leukemia (including small lymphocytic lymphoma).